PMDA approval: Arranon G Injection 250 mg
Applicant: GlaxoSmithKline K.K. Approval date: 2007-10-19. Type: New approval. Active ingredient(s): Nelarabine.
Notes: A drug containing a new active ingredient indicated for the treatment of relapsed or refractory T-cell acute lymphoblastic leukemia and T-cell lymphoblastic lymphoma. [Orphan drug]